PMDA approval: Imfinzi Injection 120 mg Imfinzi Injection 500 mg
Applicant: AstraZeneca K.K. Approval date: 2023-11-24. Type: Partial change. Active ingredient(s): Durvalumab (genetical recombination).
Notes: Drugs with a new dosage indicated for the maintenance treatment of locally-advanced, unresectable non-small cell lung cancer following definitive chemoradiation therapy.